PMDA approval: Trulicity Subcutaneous Injection 0.75 mg Ateos
Applicant: Eli Lilly Japan K.K. Approval date: 2015-07-03. Type: New approval. Active ingredient(s): Dulaglutide (genetical recombination).
Notes: A drug with a new active ingredient indicated for the treatment of type 2 diabetes mellitus.